PMDA approval: Stivarga Tablets 40 mg
Applicant: Bayer Yakuhin, Ltd. Approval date: 2013-08-20. Type: Partial change. Active ingredient(s): Regorafenib hydrate.
Notes: A drug with a new additional indication for the treatment of gastrointestinal stromal tumor which has progressed after cancer chemotherapy. [Priority review]